PMDA approval: Zytiga Tablets 250 mg
Applicant: Janssen Pharmaceutical K.K. Approval date: 2018-02-16. Type: Partial change. Active ingredient(s): Abiraterone acetate.
Notes: A drug with a new additional indication for the treatment of prostate cancer in patients with high-risk prognostic factors who have not received previous endocrine therapy. [Priority review]